PMDA approval: Oxycontin TR Tablets 5 mg Oxycontin TR Tablets10 mg Oxycontin TR Tablets 20 mg Oxycontin TR Tablets 40 mg
Applicant: Shionogi Pharma Co., Ltd. Approval date: 2020-10-29. Type: Partial change. Active ingredient(s): Oxycodone hydrochloride hydrate.
Notes: Drugs with a new indication and a new dosage for analgesia in moderate to severe chronic pain which cannot be managed by treatment with non-opioid analgesics or other opioid analgesics.